PMDA approval: Dacarbazine Injection 100
Applicant: Kyowa Hakko Kirin Co., Ltd. Approval date: 2013-03-25. Type: Partial change. Active ingredient(s): Dacarbazine.
Notes: A drug with a new additional indication and a new dosage for the treatment of pheochromocytoma. [Public knowledge-based application after PAFSC's preliminary assessment]